PMDA approval: Taxotere 20 mg for I.V. Infusion Taxotere 80 mg for I.V. Infusion
Applicant: Sanofi-Aventis K.K. Approval date: 2010-11-16. Type: Partial change. Active ingredient(s): Docetaxel hydrate.
Notes: Drugs with new dosages for the treatment of breast cancer, non-small cell lung cancer, gastric cancer, head and neck cancer and ovarian cancer.